PMDA approval: Zyprexa Tablets 2.5 mg Zyprexa Tablets 5 mg Zyprexa Tablets 10 mg Zyprexa Fine Granule 1% Zyprexa Zydis Tablets 5 mg Zyprexa Zydis Tablets 10 mg
Applicant: Eli Lilly Japan K.K. Approval date: 2012-02-22. Type: Partial change. Active ingredient(s): Olanzapine.
Notes: Drugs with a new additional indication and a new dosage for the improvement of depressive symptoms in patients with bipolar disorder.